PMDA approval: Darvias Injection 135 mg
Applicant: Solasia Pharma K.K. Approval date: 2022-06-20. Type: New approval. Active ingredient(s): Darinaparsin.
Notes: A drug with a new active ingredient indicated for the treatment of relapsed or refractory peripheral T-cell lymphoma.